PMDA approval: Rystiggo for S.C. Injection 280 mg
Applicant: UCB Japan Co., Ltd. Approval date: 2023-09-25. Type: New approval. Active ingredient(s): Rozanolixizumab (genetical recombination).
Notes: A drug with a new active ingredient indicated for the treatment of generalized myasthenia gravis (for use only in patients who have not sufficiently responded to steroids or other immunosuppressants). [Orphan drug]